PMDA approval: Samsca Tablets 7.5 mg Samsca Tablets 15 mg Samsca Tablets 30 mg
Applicant: Otsuka Pharmaceutical Co., Ltd. Approval date: 2014-03-24. Type: Partial change. Active ingredient(s): Tolvaptan.
Notes: Drugs with a new additional indication and a new dosage, and a drug with a newly-added dosage form indicated for inhibiting the progression of autosomal dominant polycystic kidney disease in patients whose kidney volume already have increased and enlarged at a rapid rate. [Orphan drug]